PMDA approval: L-Cartin Tablets 100 mg L-Cartin Tablets 300 mg
Applicant: Otsuka Pharmaceutical Co., Ltd. Approval date: 2011-03-10. Type: Partial change. Active ingredient(s): Levocarnitine chloride.
Notes: Drugs with a new additional indication and new dosages for the treatment of carnitine deficiency. [Public knowledge-based application after PAFSC's preliminary assessment]